PMDA approval: Nubeqa Tablets 300 mg
Applicant: Bayer Yakuhin, Ltd. Approval date: 2023-02-24. Type: Partial change. Active ingredient(s): Darolutamide.
Notes: A drug with a new indication and a new dosage for the treatment of metastatic prostate cancer.